PMDA approval: Relumina Tablets 40 mg
Applicant: ASKA Pharmaceutical Co., Ltd. Approval date: 2021-12-24. Type: Partial change. Active ingredient(s): Relugolix.
Notes: A drug with a new indication for the alleviation of pain associated with endometriosis.